PMDA approval: Bepio Wash Gel 5%
Applicant: Maruho Co., Ltd. Approval date: 2025-03-27. Type: New approval. Active ingredient(s): Benzoyl peroxide.
Notes: A drug with a new dosage in an additional dosage form indicated for the treatment of acne vulgaris.